PMDA approval: Teribone Inj. 56.5 μg
Applicant: Asahi Kasei Pharma Corporation. Approval date: 2011-09-26. Type: New approval. Active ingredient(s): Teriparatide acetate.
Notes: A drug with a new route of administration, a new indication, and a new dosage indicated for the treatment of osteoporosis with high risk of fracture.